PMDA approval: Palynziq Subcutaneous Injection 2.5 mg Palynziq Subcutaneous Injection 10 mg Palynziq Subcutaneous Injection 20 mg
Applicant: BioMarin Pharmaceutical Japan K.K. Approval date: 2023-03-27. Type: New approval. Active ingredient(s): Pegvaliase (genetical recombination).
Notes: Drugs with a new active ingredient indicated for the treatment of phenylketonuria. [Orphan drug]